PMDA approval: Cancidas for Intravenous Drip Infusion 50 mg Cancidas for Intravenous Drip Infusion 70 mg
Applicant: MSD K.K. Approval date: 2014-12-18. Type: Partial change. Active ingredient(s): Caspofungin acetate.
Notes: Drugs with a new additional pediatric dosage indicated for the treatment of febrile neutropenia with suspected fungal infection, esophageal candidiasis, invasive candidiasis, and aspergillosis caused by Candida or Aspergillus.